PMDA approval: Lytgobi tablets 4 mg
Applicant: Taiho Pharmaceutical Co., Ltd. Approval date: 2023-06-26. Type: New approval. Active ingredient(s): Futibatinib.
Notes: A drug with a new active ingredient indicated for the treatment of unresectable FGFR2 fusion gene-positive biliary tract cancer that has progressed after cancer chemotherapy.